PMDA approval: (1) Trelegy 200 Ellipta 30 doses (2) Trelegy 200 Ellipta 14 doses (3) Trelegy 100 Ellipta 30 doses (4) Trelegy 100 Ellipta 14 doses
Applicant: GlaxoSmithKline K.K. Approval date: 2020-11-27. Type: New approval. Active ingredient(s): Fluticasone furoate/Umeclidinium bromide/Vilanterol trifenatate.
Notes: (1), (2) New combination drugs indicated for the treatment of bronchial asthma (when a combination treatment of an inhaled steroid, a long-acting inhaled anticholinergic agent and a long-acting beta-2 agonist is needed). (3), (4) Drugs with a new indication and a new dosage for the treatment of bronchial asthma (when a combination treatment of an inhaled steroid, a long-acting inhaled anticholinergic agent and a long-acting beta-2 agonist is needed).